PMDA approval: Busulfex Injection 60 mg
Applicant: Otsuka Pharmaceutical Co., Ltd. Approval date: 2018-09-21. Type: Partial change. Active ingredient(s): Busulfan.
Notes: A drug with a new additional once-daily dosage indicated for a conditioning regimen prior to allogeneic hematopoietic stem cell transplantation, and autologous hematopoietic stem cell transplantation for Ewing's sarcoma (ES) family tumors and neuroblastoma. [Public knowledge-based application after PAFSC's preliminary assessment]